PMDA approval: Besremi Subcutaneous Injection 250 µg Syringes Besremi Subcutaneous Injection 500 µg Syringes
Applicant: PharmaEssence Japan K.K. Approval date: 2023-03-27. Type: New approval. Active ingredient(s): Ropeginterferon alfa-2b (genetical recombination).
Notes: Drugs with a new active ingredient indicated for the treatment of polycythaemia vera (use only when conventional therapies are not sufficiently effective or inappropriate).